PMDA approval: COVID-19 Vaccine Moderna Intramuscular Injection
Applicant: Takeda Pharmaceutical Company Limited. Approval date: 2021-05-21. Type: New approval. Active ingredient(s): CX-024414.
Notes: A drug with a new active ingredient indicated for the prevention of disease caused by SARS-CoV-2 infection (COVID-19). [Drug pertaining to Special Approval for Emergency]